PMDA approval: Freeze-dried Live Attenuated Measles, Rubella Combined Vaccine "Hokken"
Applicant: The Kitasato Institute. Approval date: 2011-01-14. Type: New approval. Active ingredient(s): Freeze-dried live attenuated measles, rubella combined vaccine.
Notes: A drug with a new active ingredient indicated for the prevention of measles and rubella.